PMDA approval: Avolve Capsules 0.5 mg
Applicant: GlaxoSmithKline K.K. Approval date: 2009-07-07. Type: New approval. Active ingredient(s): Dutasteride.
Notes: A drug with a new active ingredient indicated for the treatment of benign prostatic hyperplasia.